PMDA approval: Adsorbed Cell Culture-derived Influenza Vaccine H5N1 for Intramuscular Injection 30μg/mL “Kitasato Daiichi Sankyo” Adsorbed Cell Culture-derived Influenza Vaccine H5N1 for Intramuscular Injection 60μg/mL “Kitasato Daiichi sankyo”
Applicant: Kitasato Daiichi Sankyo Vaccine Co., Ltd. Approval date: 2014-03-24. Type: New approval. Active ingredient(s): Adsorbed cell culture-derived influenza vaccine (H5N1).
Notes: Drugs with a new active ingredient indicated for the prevention of pandemic influenza (H5N1). [Orphan drug]